PMDA approval: Onoact for Intravenous Infusion 50 mg Onoact for Intravenous Infusion 150 mg
Applicant: Ono Pharmaceutical Co., Ltd. Approval date: 2019-03-26. Type: Partial change. Active ingredient(s): Landiolol hydrochloride.
Notes: Drugs with new additional indications and a new dosage for the following life-threatening cardiac arrhythmia in refractory and urgent cases: ventricular fibrillation and hemodynamically unstable ventricular tachycardia. [Orphan drug]